PMDA approval: Tecentriq Intravenous Infusion 1200 mg
Applicant: Chugai Pharmaceutical Co., Ltd. Approval date: 2019-11-22. Type: Partial change. Active ingredient(s): Atezolizumab (genetical recombination).
Notes: A drug with a new dosage indicated for the treatment of unresectable advanced or recurrent non-small-cell lung cancer.